PMDA approval: Gazyva Intravenous Infusion 1000 mg
Applicant: Chugai Pharmaceutical Co., Ltd. Approval date: 2018-07-02. Type: New approval. Active ingredient(s): Obinutuzumab (genetical recombination).
Notes: A drug with a new active ingredient indicated for the treatment of CD20-positive follicular lymphoma.